PMDA approval: Seebri Inhalation Capsules 50 μg
Applicant: Novartis Pharma K.K. Approval date: 2012-09-28. Type: New approval. Active ingredient(s): Glycopyrronium bromide.
Notes: A drug with a new active ingredient indicated for the relief of symptoms secondary to airway obstructive disorder in chronic obstructive pulmonary disease (chronic bronchitis, emphysema).